PMDA approval: FDGscan Injectable
Applicant: Nihon Medi-Physics Co., Ltd. Approval date: 2018-02-16. Type: Partial change. Active ingredient(s): Fludeoxyglucose (18F).
Notes: A drug with a new additional indication for visualization of inflammatory sites in the diagnosis of large-vessel vasculitis. [Public knowledge-based application after PAFSC's preliminary assessment]